PMDA approval: Nexletol Tablets 180 mg
Applicant: Otsuka Pharmaceutical Co., Ltd. Approval date: 2025-09-19. Type: New approval. Active ingredient(s): Bempedoic acid.
Notes: A drug with a new active ingredient indicated for the treatment of hypercholesterolemia and familial hypercholesterolemia.